PMDA approval: Exforge Combination Tablets
Applicant: Novartis Pharma K.K. Approval date: 2010-01-20. Type: New approval. Active ingredient(s): Valsartan/amlodipine besylate.
Notes: A new combination drug indicated for the treatment of hypertension.